PMDA approval: Cylocide Injection 20 mg Cylocide Injection 40 mg Cylocide Injection 60 mg Cylocide Injection 100 mg Cylocide Injection 200 mg
Applicant: Nippon Shinyaku Co., Ltd. Approval date: 2014-12-18. Type: Partial change. Active ingredient(s): Cytarabine.
Notes: Drugs with a new route of administration indicated for the treatment of acute leukemia (including acute erythroid leukemia and blast crisis of chronic myelogenous leukemia). [Expedited review]